PMDA approval: Kenketsu Venilon-I for Intravenous Injection 500 mg Kenketsu Venilon-I for Intravenous Injection 1000 mg Kenketsu Venilon-I for Intravenous Injection 2500 mg Kenketsu Venilon-I for Intravenous Injection 5000 mg
Applicant: KM Biologics Co., Ltd. Approval date: 2019-08-22. Type: Partial change. Active ingredient(s): Freeze-dried sulfonated human normal immunoglobulin.
Notes: Drugs with a new additional indication and a new dosage for the improvement of muscle weakness in patients with chronic inflammatory demyelinating polyneuropathy (including multifocal motor neuropathy).